PMDA approval: Treakisym Injection 100 mg
Applicant: SymBio Pharmaceuticals Limited. Approval date: 2010-10-27. Type: New approval. Active ingredient(s): Bendamustine hydrochloride.
Notes: A drug with a new active ingredient indicated for the treatment of relapsed or refractory indolent B-cell non-Hodgkin’s lymphoma and mantle cell lymphoma. [Orphan drug]